PMDA approval: Irtra Combination Tablets LD Irtra Combination Tablets HD
Applicant: Shionogi & Co., Ltd. Approval date: 2013-06-28. Type: New approval. Active ingredient(s): Irbesartan/ trichlormethiazide.
Notes: New combination drugs indicated for the treatment of hypertension.